PMDA approval: Forteo S.C. Injection Kit 600 μg Forteo S.C. Injection Cart 600 μg
Applicant: Eli Lilly Japan K.K. Approval date: 2010-07-23. Type: New approval. Active ingredient(s): Teriparatide (genetical recombination).
Notes: Drugs with a new active ingredient indicated for the treatment of osteoporosis with a high risk of fracture.